PMDA approval: Jakavi Tablets 5 mg
Applicant: Novartis Pharma K.K. Approval date: 2014-07-04. Type: New approval. Active ingredient(s): Ruxolitinib phosphate.
Notes: A drug with a new active ingredient indicated for the treatment of myelofibrosis. [Orphan drug]